PMDA approval: Tryptanol Tablets 10 Tryptanol Tablets 25 (Nichi-Iko Pharmaceutical Co., Ltd.) Amitriptyline Hydrochloride Tablets 10 mg "Sawai" Amitriptyline Hydrochloride Tablets 25 mg "Sawai"
Applicant: Sawai Pharmaceutical Co., Ltd. Approval date: 2016-02-29. Type: Partial change. Active ingredient(s): Amitriptyline hydrochloride.
Notes: Drugs with a new additional indication and a new dosage for the treatment of peripheral neuropathic pain. [Public knowledge-based application after preliminary assessment by the Pharmaceutical Affairs and Food Sanitation Council (PAFSC)]